PMDA approval: Methapain Tablet 5 mg Methapain Tablet 10 mg
Applicant: Teikoku Seiyaku Co., Ltd. Approval date: 2012-09-28. Type: New approval. Active ingredient(s): Methadone hydrochloride.
Notes: Drugs with a new active ingredient indicated for achieving analgesia of moderate to severe pain associated with various types of cancer which can not be managed by treatment with other strong opioid analgesics.